PMDA approval: Rapiacta 300 mg Bag for Intravenous Drip Infusion Rapiacta 150 mg Vial for Intravenous Drip Infusion
Applicant: Shionogi & Co., Ltd. Approval date: 2010-10-27. Type: Partial change. Active ingredient(s): Peramivir hydrate.
Notes: Drugs with new additional pediatric dosages for influenza A or B virus infection.